PMDA approval: Ferinject Solution for Injection/Infusion 500 mg
Applicant: Zeria Pharmaceutical Co., Ltd. Approval date: 2019-03-26. Type: New approval. Active ingredient(s): Ferric carboxymaltose.
Notes: A drug with a new active ingredient indicated for the treatment of iron-deficiency anemia.